PMDA approval: Zaltrap 100 mg I.V. Infusion Zaltrap 200 mg I.V. Infusion
Applicant: Sanofi K.K. Approval date: 2017-03-30. Type: New approval. Active ingredient(s): Aflibercept beta (genetical recombination).
Notes: Drugs with a new active ingredient indicated for the treatment of unresectable advanced or recurrent colorectal cancer.